PMDA approval: Jadenu Granules Sachet 90 mg Jadenu Granules Sachet 360 mg
Applicant: Novartis Pharma K.K. Approval date: 2017-07-03. Type: New approval. Active ingredient(s): Deferasirox.
Notes: Drugs in a new dosage form indicated for the treatment of chronic iron overload due to blood transfusions (in patients for whom injection of iron chelating agents is inappropriate).